PMDA approval: Denosumab BS 120 mg RM for S.C. Injection "F"
Applicant: Fuji Pharma Co., Ltd. Approval date: 2025-09-19. Type: New approval. Active ingredient(s): Denosumab (genetical recombination) [denosumab biosimilar 1].
Notes: A biosimilar indicated for the treatment of bone lesions due to multiple myeloma and bone metastasis of solid tumor.